PMDA approval: Kaytwo Syrup 0.2%
Applicant: Sannova Co., Ltd. Approval date: 2012-05-25. Type: Partial change. Active ingredient(s): Menatetrenone.
Notes: A drug with a new additional indication and a new dosage for the prevention of vitamin K-deficiency hemorrhage in neonates/infants. [Public knowledge-based application after preliminary assessment by the Pharmaceutical Affairs and Food Sanitation Council (PAFSC)]